PMDA approval: Remitch Capsules 2.5 µg
Applicant: Toray Industries, Inc. Approval date: 2009-01-21. Type: New approval. Active ingredient(s): Nalfurafine hydrochloride.
Notes: A drug containing a new active ingredient indicated for the treatment of pruritus in hemodialysis patients (for use only when conventional therapies are not sufficiently effective).